PMDA approval: Locametz Kit
Applicant: Novartis Pharma K.K. Approval date: 2025-09-19. Type: New approval. Active ingredient(s): Gozetotide.
Notes: A drug with a new active ingredient indicated for the support of selection of patients with prostate cancer, for whom PSMA-targeted therapy is indicated.